PMDA approval: Koselugo Capsules 10 mg Koselugo Capsules 25 mg
Applicant: Alexion Pharma GK. Approval date: 2025-08-25. Type: Partial change. Active ingredient(s): Selmetinib sulfate.
Notes: Drugs with a new dosage indicated for the treatment of plexiform neurofibroma in patients with neurofibromatosis type 1. [Orphan drug]